PMDA approval: Rezurock Tablets 200 mg
Applicant: Meiji Seika Pharma Co., Ltd. Approval date: 2024-03-26. Type: New approval. Active ingredient(s): Belumosudil mesilate.
Notes: A drug with a new active ingredient indicated for the treatment of chronic graft-versus-host disease after hematopoietic stem cell transplantation (for use when steroid drugs are not sufficiently effective). [Orphan drug]